PMDA approval: Adsorbed Pandemic Influenza Vaccine (H5N1) "Biken"
Applicant: The Research Foundation for Microbial Diseases of Osaka University. Approval date: 2007-10-19. Type: New approval. Active ingredient(s): Inactivated influenza virus, H5N1 (whole virion).
Notes: A drug containing a new active ingredient indicated for the prevention of pandemic influenza (H5N1). [Orphan drug]